PMDA approval: Talion Tablets 5 mg Talion Tablets 10 mg Talion OD Tablets 5 mg Talion OD Tablets 10 mg
Applicant: Mitsubishi Tanabe Pharma Corporation. Approval date: 2015-05-26. Type: Partial change. Active ingredient(s): Bepotastine besilate.
Notes: Drugs with a new additional pediatric dosage indicated for the treatment of allergic rhinitis, urticaria, and itching associated with skin diseases (eczema/dermatitis, cutaneous pruritus).